PMDA approval: Streptomycin Sulfate 1 g ”Meiji” for Injection
Applicant: Meiji Seika Pharma Co., Ltd. Approval date: 2014-02-21. Type: Partial change. Active ingredient(s): Streptomycin sulfate.
Notes: A drug with a new additional indication and new dosage for the treatment of non-tuberculous mycobacterial infection including mycobacterium avium complex (MAC) caused by streptomycin-sensitive mycobacterium as its applicable microorganism. [Public knowledge-based application after PAFSC's preliminary assessment]